PMDA approval: Metact Combination Tablets LD Metact Combination Tablets HD
Applicant: Takeda Pharmaceutical Company Limited. Approval date: 2010-04-16. Type: New approval. Active ingredient(s): Pioglitazone hydrochloride/metformin hydrochloride.
Notes: New combination drugs indicated for the treatment of type 2 diabetes (only when a concomitant use of pioglitazone hydrochloride with metformin hydrochloride is deemed appropriate).